PMDA approval: Lucentis solution for intravitreal injection 2.3 mg/0.23 mL
Applicant: Novartis Pharma K.K. Approval date: 2014-02-21. Type: Partial change. Active ingredient(s): Ranibizumab (genetical recombination).
Notes: A drug with a new additional indication for the treatment of diabetic macular edema.